PMDA approval: Regpara Tablets 25 mg Regpara Tablets 75 mg
Applicant: Kyowa Hakko Kirin Co., Ltd. Approval date: 2014-02-21. Type: Partial change. Active ingredient(s): Cinacalcet hydrochloride.
Notes: Drugs with a new additional indication and a new dosage for the treatment of hypercalcemia in patients with parathyroid carcinoma, and hypercalcemia in patients with primary hyperparathyroidism (HPT) who are unable to undergo parathyroidectomy or who experience recurrent primary HPT after the surgery. [Orphan drug]